PMDA approval: NovoRapid 50 Mix Penfill NovoRapid 50 Mix FlexPen
Applicant: Novo Nordisk Pharma Ltd. Approval date: 2009-09-18. Type: New approval. Active ingredient(s): Insulin aspart (genetical recombination).
Notes: Drugs with a new dosage and in an additional dosage form indicated for the treatment of diabetes mellitus where insulin therapy is indicated.